PMDA approval: Jaypirca Tablets 50 mg Jaypirca Tablets 100 mg
Applicant: Eli Lilly Japan K.K. Approval date: 2025-09-19. Type: Partial change. Active ingredient(s): Pirtobrutinib.
Notes: Drugs with a new indication for the treatment of relapsed or refractory chronic lymphocytic leukemia (including small lymphocytic lymphoma) with resistant or intolerant to other BTK inhibitors.